PMDA approval: Libtayo I.V. Infusion 350 mg
Applicant: Sanofi K.K. Approval date: 2022-12-23. Type: New approval. Active ingredient(s): Cemiplimab (genetical recombination).
Notes: A drug with a new active ingredient indicated for the treatment of advanced or recurrent cervical cancer that has progressed after cancer chemotherapy.